PMDA approval: Warfarin Tablets 0.5 mg Warfarin Tablets 1 mg Warfarin Tablets 5 mg
Applicant: Eisai Co., Ltd. Approval date: 2011-02-23. Type: Partial change. Active ingredient(s): Warfarin potassium.
Notes: Drugs with new additional pediatric dosages. These drugs are indicated for the treatment and prophylaxis of thromboembolism (e.g., phlebothrombosis, myocardial infarction, pulmonary embolism, brain embolism, slowly-progressive cerebral thrombosis). [Public knowledge-based application after PAFSC's preliminary assessment]